PMDA approval: Adzynma Intravenous 1500
Applicant: Takeda Pharmaceutical Company Limited. Approval date: 2024-03-26. Type: New approval. Active ingredient(s): Apadamtase alfa (genetical recombination)/ Cinaxadamtase alfa (genetical recombination).
Notes: A drug with a new active ingredient indicated for the treatment of congenital thrombotic thrombocytopenic purpura. [Orphan drug]